PMDA approval: Riamet Combination Tablets
Applicant: Novartis Pharma K.K. Approval date: 2016-12-19. Type: New approval. Active ingredient(s): Artemether/ Lumefantrine.
Notes: A new combination drug with new active ingredients indicated for the treatment of malaria.